PMDA approval: Xolair for S.C. Injection 150 mg Xolair for S.C. Injection 75 mg
Applicant: Novartis Pharma K.K. Approval date: 2017-03-24. Type: Partial change. Active ingredient(s): Omalizumab (genetical recombination).
Notes: Drugs with a new additional indication and a new dosage for the treatment of chronic idiopathic urticaria (for use only in patients who have not responded sufficiently to conventional therapies).